PMDA approval: Ozex Fine Granules 15% for Pediatric
Applicant: Toyama Chemical Co., Ltd. Approval date: 2009-10-16. Type: New approval. Active ingredient(s): Tosufloxacin tosilate hydrate.
Notes: A drug with new additional indications and a new dosage and in a new dosage form for the treatment of pneumonia, cholera, otitis media, and anthrax in children.